PMDA approval: Lenvima Capsules 4 mg Lenvima Capsules 10 mg
Applicant: Eisai Co., Ltd. Approval date: 2022-02-25. Type: Partial change. Active ingredient(s): Lenvatinib mesilate.
Notes: Drugs with a new indication and a new dosage for the treatment of unresectable or metastatic renal cell carcinoma.